PMDA approval: Kenketu Glovenin-I for I.V. Injection 2500 mg Kenketu Glovenin-I for I.V. Injection 500 mg Kenketu Glovenin-I for I.V. Injection 5000 mg
Applicant: Nihon Pharmaceutical Co., Ltd. Approval date: 2016-12-19. Type: Partial change. Active ingredient(s): Polyethylene glycol treated human normal immunoglobulin G.
Notes: Drugs with a new additional indication and a new dosage for inhibiting progression of motor disability due to chronic inflammatory demyelinating polyneuropathy (including multifocal motor neuropathy) (in the cases where patients show an improvement in their acute phase treatment) [Orphan drug]